PMDA approval: Neutrogin for Injection 50 μg Neutrogin for Injection 100 μg Neutrogin for Injection 250 μg
Applicant: Chugai Pharmaceutical Co., Ltd. Approval date: 2022-06-20. Type: Partial change. Active ingredient(s): Lenograstim (genetical recombination).
Notes: Drugs with a new indication and a new dosage for use as an adjunctive therapy with antineoplastic agents for the treatment of relapsed or refractory acute myeloid leukemia. [Public knowledge-based application after PAFSC's preliminary assessment]